PMDA approval: Enhertu for Intravenous Drip Infusion 100 mg
Applicant: Daiichi Sankyo Company, Limited. Approval date: 2022-11-24. Type: Partial change. Active ingredient(s): Trastuzumab deruxtecan (genetical recombination).
Notes: A drug with a new indication for the treatment of unresectable or recurrent HER2-positive breast cancer in patients who have previously been treated with chemotherapy.